PMDA approval: Tarceva Tablet 25 mg Tarceva Tablet 100 mg Tarceva Tablet 150 mg
Applicant: Chugai Pharmaceutical Co., Ltd. Approval date: 2013-06-14. Type: Partial change. Active ingredient(s): Erlotinib hydrochloride.
Notes: Drugs with a new additional indication and a new dosage for the treatment of unresectable advanced or recurrent non-small cell lung cancer with EGFR gene mutation in patients who have not been treated with chemotherapy.